PMDA approval: Zosyn for Intravenous Injection 2.25 Zosyn for Intravenous Injection 4.5 Zosyn for I.V. Infusion bag 4.5
Applicant: Taiho Pharmaceutical Co., Ltd. Approval date: 2015-06-26. Type: Partial change. Active ingredient(s): Tazobactam/ Piperacillin hydrate.
Notes: Drugs with a new additional indication and a new dosage for the treatment of febrile neutropenia.